PMDA approval: (1) Optiray 350 Injection 20 mL (2) Optiray 350 Injection 50 mL (3) Optiray 350 Injection 100 mL (4) Optiray 350 Injection Syringe 100 mL (5) Optiray 350 Injection Syringe 135 mL
Applicant: Fuji Pharma Co., Ltd. Approval date: 2017-03-30. Type: Partial change. Active ingredient(s): Ioversol.
Notes: Drugs with a new additional indication and a new dosage, and an additional dosage form used as contrast media in abdominal computed tomography.